PMDA approval: Omepral Tablets 10 (AstraZeneca K.K.) Omeprazon Tablets 10 mg
Applicant: Mitsubishi Pharma Corporation. Approval date: 2007-05-24. Type: Partial change. Active ingredient(s): Omeprazole.
Notes: Drugs with a new indication and dosage for the treatment of non-erosive reflux disease.